PMDA approval: DuoTrav Combination Ophthalmic Solution
Applicant: Alcon Japan Ltd. Approval date: 2010-04-16. Type: New approval. Active ingredient(s): Travoprost/timolol maleate.
Notes: A new combination drug indicated for the treatment of glaucoma and ocular hypertension.